PMDA approval: (1) Fasenra Subcutaneous Injection 30 mg Syringe (2) Fasenra Subcutaneous Injection 30 mg Pen
Applicant: AstraZeneca K.K. Approval date: 2024-12-27. Type: Partial change. Active ingredient(s): Benralizumab (genetical recombination).
Notes: (1) A drug with a new indication and a new dosage and (2) a drug with a new indication and a new dosage in an additional dosage form, indicated for the treatment of eosinophilic granulomatosis with polyangiitis in patients who have not responded sufficiently to conventional therapies.